PMDA approval: Anerem 50 mg for I.V. Injection
Applicant: Mundipharma K.K. Approval date: 2020-01-23. Type: New approval. Active ingredient(s): Remimazolam besilate.
Notes: A drug with a new active ingredient indicated for the induction and maintenance of general anesthesia.